PMDA approval: Moizerto Ointment 0.3% Moizerto Ointment 1%
Applicant: Otsuka Pharmaceutical Co., Ltd. Approval date: 2021-09-27. Type: New approval. Active ingredient(s): Difamilast.
Notes: Drugs with a new active ingredient indicated for the treatment of atopic dermatitis.